PMDA approval: Aibeta Combination Ophthalmic Solution
Applicant: Senju Pharmaceutical Co., Ltd. Approval date: 2019-09-20. Type: New approval. Active ingredient(s): Brimonidine tartrate/Timolol maleate.
Notes: A new combination drug indicated for the treatment of glaucoma and ocular hypertension in patients who have not responded sufficiently to other anti-glaucoma drugs.